PMDA approval: Amvuttra Subcutaneous Injection 25 mg Syringe
Applicant: Alnylam Japan, K.K. Approval date: 2022-09-26. Type: New approval. Active ingredient(s): Vutrisiran sodium.
Notes: A drug with a new active ingredient indicated for the treatment of transthyretin familial amyloid polyneuropathy. [Orphan drug]